PMDA approval: Enerzair Inhalation Capsules Medium Dose Enerzair Inhalation Capsules High Dose
Applicant: Novartis Pharma K.K. Approval date: 2020-06-29. Type: New approval. Active ingredient(s): Indacaterol acetate/Glycopyrronium bromide/Mometasone furoate.
Notes: New combination drugs with a new active ingredient indicated for the treatment of bronchial asthma (when a combination treatment of an inhaled steroid, a long-acting beta-2 agonist and a long-acting inhaled anticholinergic agent is needed).